PMDA approval: Rinvoq Tablets 7.5 mg Rinvoq Tablets 15 mg Rinvoq Tablets 30 mg
Applicant: AbbVie GK. Approval date: 2024-09-24. Type: Partial change. Active ingredient(s): Upadacitinib hydrate.
Notes: Drugs with a new additional pediatric dosage indicated for the treatment of atopic dermatitis in patients who have not responded sufficiently to conventional treatments.